PMDA approval: Keytruda Injection 20 mg Keytruda Injection 100 mg
Applicant: MSD K.K. Approval date: 2016-09-28. Type: New approval. Active ingredient(s): Pembrolizumab (genetical recombination).
Notes: Drugs with a new active ingredient indicated for the treatment of unresectable melanoma. [Orphan drug]